PMDA approval: Gran Injection 75 Gran Injection 150 Gran Injection M300 Gran Syringe 75 Gran Syringe 150 Gran Syringe M300
Applicant: Kyowa Kirin Co., Ltd. Approval date: 2021-06-23. Type: Partial change. Active ingredient(s): Filgrastim (genetical recombination).
Notes: Drugs with a new indication and a new dosage for the enhancement of an antitumor effect of dinutuximab (genetical recombination) for neuroblastoma. [Expedited review]